PMDA approval: Tecentriq Intravenous Infusion 1200 mg
Applicant: Chugai Pharmaceutical Co., Ltd. Approval date: 2020-09-25. Type: Partial change. Active ingredient(s): Atezolizumab (genetical recombination).
Notes: A drug with a new indication and a new dosage for the treatment of unresectable hepatocellular carcinoma. [Priority review]